PMDA approval: Solu-Medrol for Intravenous Use 40 mg Solu-Medrol for Intravenous Use 125 mg Solu-Medrol for Intravenous Use 500 mg Solu-Medrol for Intravenous Use 1000 mg (Pfizer Japan Inc.) Sol-Melcort for Injection 40 Sol-Melcort for Injection 125 Sol-Melcort for Injection 500 Sol-Melcort for Injection 1,000
Applicant: Fuji Pharma Co., Ltd. Approval date: 2011-05-20. Type: Partial change. Active ingredient(s): Methylprednisolone sodium succinate.
Notes: Drugs with a new additional indication and a new dosage for the treatment of nephrotic syndrome. [Public knowledge-based application after preliminary assessment by the Pharmaceutical Affairs and Food Sanitation Council (PAFSC)]